PMDA approval: Emla Cream
Applicant: Sato Pharmaceutical Co., Ltd. Approval date: 2015-06-26. Type: Partial change. Active ingredient(s): Lidocaine/Propitocain e.
Notes: A drug with a new additional indication and a new dosage for adults indicated for the relief of pain during pricking with injection needles and intravenous indwelling needles, and with a new pediatric dosage indicated for the relief of pain during skin laser radiation therapy and pricking with injection needles and intravenous indwelling needles.